PMDA approval: Herceptin Injection 60 Herceptin Injection 150
Applicant: Chugai Pharmaceutical Co., Ltd. Approval date: 2008-02-29. Type: Partial change. Active ingredient(s): Trastuzumab (genetical recombination).
Notes: Drugs with a new indication and dosage for use as a postoperative adjuvant chemotherapy in patients with HER2-overexpressing breast cancer. [Priority review]